PMDA approval: Trerief Tablets 25 mg
Applicant: Dainippon Sumitomo Pharma Co., Ltd. Approval date: 2009-01-21. Type: New approval. Active ingredient(s): Zonisamide.
Notes: A drug with a new indication and a new dosage for the treatment of Parkinson's disease (in patients who have been treated with other anti-Parkinson drugs in combination with a levodopa-containing agent, but sufficient therapeutic effect was not obtained).